PMDA approval: Rotarix Oral Solution
Applicant: GlaxoSmithKline K.K. Approval date: 2011-07-01. Type: New approval. Active ingredient(s): Live attenuated human rotavirus vaccine, oral.
Notes: A drug with a new active ingredient indicated for the prevention of gastroenteritis due to rotavirus.